PMDA approval: Dysval Capsules 40 mg
Applicant: Mitsubishi Tanabe Pharma Corporation. Approval date: 2022-03-28. Type: New approval. Active ingredient(s): Valbenazine tosilate.
Notes: A drug with a new active ingredient indicated for the treatment of tardive dyskinesia.